PMDA approval: Glufast Tab. 5 mg Glufast Tab. 10 mg
Applicant: Kissei Pharmaceutical Co., Ltd. Approval date: 2013-09-13. Type: Partial change. Active ingredient(s): Mitiglinide calcium hydrate.
Notes: Drugs with a new indication for the treatment of type 2 diabetes mellitus.